PMDA approval: Imuran Tablets 50 mg (GlaxoSmithKline K.K.) Azanin Tablets 50 mg
Applicant: Mitsubishi Tanabe Pharma Corporation. Approval date: 2011-05-20. Type: Partial change. Active ingredient(s): Azathioprine.
Notes: Drugs with new additional indications and a new dosage for the treatment of the following treatment-resistant rheumatic diseases: systemic vasculitis (e.g., microscopic polyangiitis, Wegener granulomatosis, polyarteritis nodosa, Churg-Strauss syndrome, aortitis syndrome), systemic lupus erythematosus (SLE), polymyositis, dermatomyositis, pachyderma, mixed connective tissue disease and refractory rheumatic disease. [Public knowledge-based application after PAFSC's preliminary assessment]